PMDA approval: Edirol Capsule 0.5 μg Edirol Capsule 0.75 μg
Applicant: Chugai Pharmaceutical Co., Ltd. Approval date: 2011-01-21. Type: New approval. Active ingredient(s): Eldecalcitol.
Notes: Drugs with a new active ingredient indicated for the treatment of osteoporosis.